PMDA approval: Concerta Tablets 36 mg
Applicant: Janssen Pharmaceutical K.K. Approval date: 2014-01-17. Type: New approval. Active ingredient(s): Methylphenidate hydrochloride.
Notes: A drug with a new indication and a new dosage in an additional dosage form indicated for the treatment of attention deficit/hyperactivity disorder (AD/HD) in adults.